PMDA approval: Kanuma Intravenous Infusion 20 mg
Applicant: Alexion Pharma G.K. Approval date: 2016-03-28. Type: New approval. Active ingredient(s): Sebelipase alfa (genetical recombination).
Notes: A drug with a new active ingredient indicated for the treatment of lysosomal acid lipase deficiency. [Orphan drug]